PMDA approval: Filgrastim BS Injection 75 μg Syringe "Mochida" Filgrastim BS Injection 150 μg Syringe "Mochida" Filgrastim BS Injection 300 μg Syringe "Mochida" (Mochida Pharmaceutical Co., Ltd.) Filgrastim BS Injection 75 μg Syringe "F" Filgrastim BS Injection 150 μg Syringe "F" Filgrastim BS Injection 300 μg Syringe "F"
Applicant: Fuji Pharma Co., Ltd. Approval date: 2012-11-21. Type: New approval. Active ingredient(s): Filgrastim (genetical recombination).
Notes: Follow-on biologics indicated for mobilization of hematopoietic stem cells to peripheral blood, promotion of increases in neutrophil count at the time of hematopoietic stem cell transplantation, and the treatment of neutropenia caused by cancer chemotherapy, neutropenia which affects the treatment of human immunodeficiency virus (HIV) infection, neutropenia associated with myelodysplastic syndrome, neutropenia associated with aplastic anemia, and congenital/idiopathic neutropenia.